PMDA approval: Hizentra 20% S.C. Injection 1 g/5 mL Hizentra 20% S.C. Injection 2 g/10 mL Hizentra 20% S.C. Injection 4 g/20 mL
Applicant: CSL Behring K.K. Approval date: 2013-09-27. Type: New approval. Active ingredient(s): pH4-treated normal human immunoglobulin (subcutaneous injection).
Notes: Drugs with a new active ingredient indicated for the treatment of agammaglobulinemia or hypogammaglobulinemia.